PMDA approval: Defitelio Injection 200 mg
Applicant: Nippon Shinyaku Co., Ltd. Approval date: 2019-06-18. Type: New approval. Active ingredient(s): Defibrotide sodium.
Notes: A drug with a new active ingredient indicated for the treatment of sinusoidal obstruction syndrome (SOS) /hepatic veno-occlusive disease (VOD). [Orphan drug]